PMDA approval: Cedartolen Sublingual Drop-Japanese Cedar Polllen 200 JAU/mL bottle Cedartolen Sublingual Drop-Japanese Cedar Polllen 2,000 JAU/mL bottle Cedartolen Sublingual Drop-Japanese Cedar Polllen 2,000 JAU/mL pack
Applicant: Torii Pharmaceutical Co., Ltd. Approval date: 2014-01-17. Type: New approval. Active ingredient(s): Standardized Japanese cedar pollen extract original solution 10,000 JAU/mL.
Notes: Drugs with a new route of administration indicated for the treatment of Japanese cedar pollinosis. (Allergen immunotherapy).